PMDA approval: Tobi Inhalation Solution 300 mg
Applicant: Novartis Pharma K.K. Approval date: 2012-09-28. Type: New approval. Active ingredient(s): Tobramycin.
Notes: A drug with a new route of administration indicated for the improvement of symptoms associated with respiratory infection caused by Pseudomonas aeruginosa in cystic fibrosis patients.